PMDA approval: Allegra 5% Dry Syrup
Applicant: Sanofi K.K. Approval date: 2014-01-17. Type: New approval. Active ingredient(s): Fexofenadine hydrochloride.
Notes: A drug with a new additional pediatric (6 months of age and older and less than 7 years of age) dosage in an additional dosage form of dry syrup. This drug is indicated for the treatment of allergic rhinitis, urticaria, and itching associated with skin diseases (eczema/dermatitis, pruritus cutaneous, atopic dermatitis).